PMDA approval: Humira 40 mg for S.C. Injection Syringe 0.4 mL Humira 80 mg for S.C. Injection Syringe 0.8 mL Humira 40 mg for S.C. Injection in pre-filled pen 0.4 mL Humira 80 mg for S.C. Injection in pre-filled pen 0.8 mL Humira 20 mg for S.C. Injection Syringe 0.2 mL
Applicant: AbbVie GK. Approval date: 2021-09-27. Type: Partial change. Active ingredient(s): Adalimumab (genetical recombination).
Notes: Drugs with a new indication and a new dosage, including a new additional pediatric dosage, for the treatment of moderate or severe ulcerative colitis (for use only in patients who have not responded sufficiently to conventional treatments).